PMDA approval: Enhertu for Intravenous Drip Infusion 100 mg
Applicant: Daiichi Sankyo Company, Limited. Approval date: 2025-08-25. Type: Partial change. Active ingredient(s): Trastuzumab deruxtecan (genetical recombination).
Notes: A drug with a new indication for the treatment of hormone receptor-positive and human epidermal growth factor receptor 2 (HER2)-low or -ultralow inoperable or recurrent breast cancer.